PMDA approval: Besponsa Injection 1 mg
Applicant: Pfizer Japan Inc. Approval date: 2018-01-19. Type: New approval. Active ingredient(s): Inotuzumab ozogamicin (genetical recombination).
Notes: A drug with a new active ingredient indicated for the treatment of relapsed or refractory CD22-positive acute lymphoblastic leukemia. [Orphan drug]